PMDA approval: Urece Tablets 0.5 mg Urece Tablets 1 mg Urece Tablets 2 mg
Applicant: Fuji Yakuhin Co., Ltd. Approval date: 2020-01-23. Type: New approval. Active ingredient(s): Dotinurad.
Notes: Drugs with a new active ingredient indicated for the treatment of gout and hyperuricemia.